PMDA approval: Miripla for Intra-arterial Injection 70 mg
Applicant: Dainippon Sumitomo Pharma Co., Ltd. Approval date: 2009-10-16. Type: New approval. Active ingredient(s): Miriplatin hydrate.
Notes: A drug with a new active ingredient indicated for lipiodolization in hepatocellular carcinoma.